PMDA approval: Mounjaro Subcutaneous Injection 2.5 mg Ateos Mounjaro Subcutaneous Injection 5 mg Ateos Mounjaro Subcutaneous Injection 7.5 mg Ateos Mounjaro Subcutaneous Injection 10 mg Ateos Mounjaro Subcutaneous Injection 12.5 mg Ateos Mounjaro Subcutaneous Injection 15 mg Ateos
Applicant: Eli Lilly Japan K.K. Approval date: 2022-09-26. Type: New approval. Active ingredient(s): Tirzepatide.
Notes: Drugs with a new active ingredient indicated for the treatment of type 2 diabetes mellitus.